PMDA approval: Brevibloc Injection 100 mg
Applicant: Maruishi Pharmaceutical Co., Ltd. Approval date: 2008-03-19. Type: Partial change. Active ingredient(s): Esmolol hydrochloride.
Notes: A drug with a new dosage indicated for the emergency treatment of supraventricular tachyarrhythmia occurring during surgery.